PMDA approval: Enbrel 10 mg for S.C. Injection Enbrel 25 mg Syringe 0.5 mL for S.C. Injection
Applicant: Wyeth K.K. Approval date: 2010-02-05. Type: Partial change. Active ingredient(s): Etanercept (genetical recombination).
Notes: Drugs with a new dosage indicated for the treatment of rheumatoid arthritis (for use only in patients who have not sufficiently responded to conventional treatments). 59 Enbrel 25 mg for S.C. Injection Enbrel 50 mg Syringe 1.0 mL for S.C. Injection (Wyeth K.K.) Change Approval